PMDA approval: Ofev Capsules 100 mg Ofev Capsules 150 mg
Applicant: Nippon Boehringer Ingelheim Co., Ltd. Approval date: 2020-05-29. Type: Partial change. Active ingredient(s): Nintedanib ethanesulfonate.
Notes: Drugs with a new indication for the treatment of chronic fibrosing interstitial lung diseases with a progressive phenotype. [Priority review]